PMDA approval: Rupafin Tablets 10 mg
Applicant: Teikoku Seiyaku Co., Ltd. Approval date: 2017-09-27. Type: New approval. Active ingredient(s): Rupatadine fumarate.
Notes: A drug with a new active ingredient indicated for the treatment of allergic rhinitis, urticaria, and itching associated with skin diseases (eczema/dermatitis, cutaneous pruritus).